PMDA approval: Erleada Tablets 60 mg
Applicant: Janssen Pharmaceutical K.K. Approval date: 2019-03-26. Type: New approval. Active ingredient(s): Apalutamide.
Notes: A drug with a new active ingredient indicated for the treatment of non-metastatic, castration-resistant prostate cancer.